PMDA approval: Lyrica Capsules 25 mg Lyrica Capsules 75 mg Lyrica Capsules 150 mg
Applicant: Pfizer Japan Inc. Approval date: 2010-04-16. Type: New approval. Active ingredient(s): Pregabalin.
Notes: Drugs with a new active ingredient indicated for the treatment of postherpetic neuralgia.